PMDA approval: Tepezza for Intravenous Infusion 500 mg
Applicant: Amgen K.K. Approval date: 2024-09-24. Type: New approval. Active ingredient(s): Teprotumumab (genetical recombination).
Notes: A drug with a new active ingredient indicated for the treatment of active thyroid eye disease. [Orphan drug]